PMDA approval: Tagrisso Tablets 40 mg Tagrisso Tablets 80 mg
Applicant: AstraZeneca K.K. Approval date: 2018-08-21. Type: Partial change. Active ingredient(s): Osimertinib mesilate.
Notes: Drugs with a new indication for the treatment of inoperable or recurrent epidermal growth factor receptor (EGFR) mutation-positive non-small cell lung cancer. [Priority review]